PMDA approval: Kostaive Intramuscular Injection
Applicant: Meiji Seika Pharma Co., Ltd. Approval date: 2023-11-28. Type: New approval. Active ingredient(s): Zapomeran.
Notes: A drug with a new active ingredient indicated for the prevention of disease caused by SARS-CoV-2 infection (COVID-19). [Priority review]